PMDA approval: Erbitux Injection 100 mg
Applicant: Merck Biopharma Co., Ltd. Approval date: 2019-09-20. Type: Partial change. Active ingredient(s): Cetuximab (genetical recombination).
Notes: A drug with a new indication for the treatment of unresectable, advanced or recurrent colon or rectal cancer with wild-type RAS.